PMDA approval: Aflibercept BS Solution 40 mg/mL for IVT Injection "NIT" Aflibercept BS Kit 40 mg/mL for IVT Injection "NIT"
Applicant: Fuji Pharma Co., Ltd. Approval date: 2025-09-19. Type: New approval. Active ingredient(s): Aflibercept (genetical recombination) [aflibercept biosimilar 2].
Notes: Biosimilars indicated for the treatment of age-related macular degeneration associated with subfoveal choroidal neovascularization, macular edema following retinal vein occlusion, and choroidal neovascularization in patients with pathologic myopia.